PMDA approval: Abraxane I.V. Infusion 100 mg
Applicant: Taiho Pharmaceutical Co., Ltd. Approval date: 2013-02-21. Type: Partial change. Active ingredient(s): Paclitaxel.
Notes: A drug with new additional indications and a new dosage for the treatment of gastric cancer and non-small cell lung cancer.